PMDA approval: NovoRapid 70 Mix Penfill NovoRapid 70 Mix FlexPen
Applicant: Novo Nordisk Pharma Ltd. Approval date: 2009-08-20. Type: New approval. Active ingredient(s): Insulin aspart (genetical recombination).
Notes: Drugs with a new dosage indicated for the treatment of diabetes mellitus where insulin therapy is indicated.